PMDA approval: Fibrinogen HT I.V. 1 g “JB”
Applicant: Japan Blood Products Organization. Approval date: 2022-03-28. Type: Partial change. Active ingredient(s): Freeze-dried human fibrinogen.
Notes: A drug with a new indication and a new dosage for the replacement of fibrinogen in patients with acquired hypofibrinogenemia associated with critical obstetrical hemorrhage. [Public knowledge-based application after PAFSC's preliminary assessment]